PMDA approval: Leuplin Pro for Injection Kit 22.5 mg
Applicant: Takeda Pharmaceutical Company Limited. Approval date: 2015-09-28. Type: New approval. Active ingredient(s): Leuprorelin acetate.
Notes: A drug in a new dosage form indicated for the treatment of prostate cancer and premenopausal breast cancer.